PMDA approval: (1) Reguneal HCa 1.5 Peritoneal Dialysis Solution Reguneal HCa 2.5 Peritoneal Dialysis Solution Reguneal HCa 4.25 Peritoneal Dialysis Solution (2) Reguneal LCa 1.5 Peritoneal Dialysis Solution Reguneal LCa 2.5 Peritoneal Dialysis Solution Reguneal LCa 4.25 Peritoneal Dialysis Solution
Applicant: Baxter Limited. Approval date: 2013-09-20. Type: New approval. Active ingredient(s): N/A for this combination drug.
Notes: Combination prescription drugs with similar formulations indicated for the treatment of (1) peritoneal dialysis in patients with chronic renal failure (used when the peritoneal dialysis is not sufficiently effective to improve hypermagnesemia), (2) peritoneal dialysis in patients with chronic renal failure (used when the peritoneal dialysis is not sufficiently effective to improve hypermagnesemia and when hypercalcaemia may occur due to the treatment with calcium preparation or active vitamin D preparation).